PMDA approval: Pivlaz I.V. Infusion liquid 150 mg
Applicant: Idorsia Pharmaceuticals Japan Ltd. Approval date: 2022-01-20. Type: New approval. Active ingredient(s): Clazosentan sodium.
Notes: A drug with a new active ingredient indicated for the prevention of cerebrovascular spasm after surgery for subarachnoid hemorrhage caused by cerebral aneurysm, and cerebral infarctions and cerebral ischemic symptoms accompanying the cerebrovascular spasm.